PMDA approval: Adcetris for Intravenous Drip Infusion 50 mg
Applicant: Takeda Pharmaceutical Company Limited. Approval date: 2018-09-21. Type: Partial change. Active ingredient(s): Brentuximab vedotin (genetical recombination).
Notes: A drug with a new indication and a new dosage for the treatment of CD30-positive Hodgkin’s lymphoma [Orphan drug]